PMDA approval: Rinvoq Tablets 7.5 mg Rinvoq Tablets 15 mg
Applicant: AbbVie GK. Approval date: 2021-05-27. Type: Partial change. Active ingredient(s): Upadacitinib hydrate.
Notes: Drugs with a new indication and a new dosage for the treatment of psoriatic arthritis in patients who have not responded sufficiently to conventional therapies.